PMDA approval: Ibrance 25 mg Tablets Ibrance 125 mg Tablets
Applicant: Pfizer Japan Inc. Approval date: 2020-01-23. Type: New approval. Active ingredient(s): Palbociclib.
Notes: Drugs with a new indication in a new dosage form for the treatment of hormone receptor (HR)-positive and human epidermal growth factor receptor 2 (HER2)-negative inoperable or recurrent breast cancer.